PMDA approval: Capvaxive for Intramuscular Injection Syringes
Applicant: MSD K.K. Approval date: 2025-08-08. Type: New approval. Active ingredient(s): Pneumococcal 21-valent conjugate vaccine (mutated diphtheria CRM197 conjugate).
Notes: A drug with a new active ingredient indicated for the prophylaxis of pneumococcal disease in the elderly or in adults who are considered to be at a high risk of pneumococcal disease.